PMDA approval: Quattrovac Subcutaneous Injection Syringe
Applicant: Kaketsuken [The Chemo-Sero-Therapeutic Research Institute]. Approval date: 2012-07-27. Type: New approval. Active ingredient(s): Adsorbed diphtheria-purified pertussis-tetanus-inactivated polio (Sabin strain) combined vaccine.
Notes: A drug with a new active ingredient indicated for the prevention of pertussis, diphtheria, tetanus, and acute poliomyelitis.